PMDA approval: Talvey Subcutaneous Injection 3 mg Talvey Subcutaneous Injection 40 mg
Applicant: Janssen Pharmaceutical K.K. Approval date: 2025-06-24. Type: New approval. Active ingredient(s): Talquetamab (genetical recombination).
Notes: Drugs with a new active ingredient indicated for the treatment of relapsed or refractory multiple myeloma (for use only if refractory or intolerant to standard therapies). [Orphan drug]